PMDA approval: Xolair for S.C. Injection
Applicant: Novartis Pharma K.K. Approval date: 2009-01-21. Type: New approval. Active ingredient(s): Omalizumab (genetical recombination).
Notes: A drug containing a new active ingredient indicated for the treatment of bronchial asthma (for use only in patients with intractable bronchial asthma whose asthmatic responses are uncontrollable with conventional therapies).